PMDA approval: Retevmo Capsules 40 mg Retevmo Capsules 80 mg
Applicant: Eli Lilly Japan K.K. Approval date: 2022-02-25. Type: Partial change. Active ingredient(s): Selpercatinib.
Notes: Drugs with a new indication and a new dosage for the treatment of RET fusion gene-positive unresectable thyroid cancer and RET gene mutation-positive unresectable medullary thyroid cancer. [Orphan drug]